PMDA approval: Tremfya Subcutaneous Injection 100 mg Syringe
Applicant: Janssen Pharmaceutical K.K. Approval date: 2018-11-21. Type: Partial change. Active ingredient(s): Guselkumab (genetical recombination).
Notes: A drug with a new additional indication for the treatment of palmoplantar pustulosis in patients who have not responded sufficiently to conventional therapies.